PMDA approval: Paxlovidpack
Applicant: Pfizer Japan Inc. Approval date: 2022-02-10. Type: New approval. Active ingredient(s): Nirmatrelvir/Ritonavir.
Notes: A drug with new active ingredients indicated for the treatment of disease caused by SARS-CoV-2 infection (COVID-19). [Drug pertaining to Special Approval for Emergency]